PMDA approval: Keytruda Injection 100 mg
Applicant: MSD K.K. Approval date: 2024-11-22. Type: Partial change. Active ingredient(s): Pembrolizumab (genetical recombination).
Notes: A drug with a new indication and a new dosage for the treatment of locally advanced cervical cancer. [Orphan drug]